PMDA approval: Allermist 27.5 μg 56 metered Nasal Spray
Applicant: GlaxoSmithKline K.K. Approval date: 2014-03-17. Type: Partial change. Active ingredient(s): Fluticasone furoate.
Notes: A drug with a new additional pediatric dosage indicated for the treatment of allergic rhinitis.